PMDA approval: Samtasu for I.V. Infusion 8 mg Samtasu for I.V. Infusion 16 mg
Applicant: Otsuka Pharmaceutical Co., Ltd. Approval date: 2022-03-28. Type: New approval. Active ingredient(s): Tolvaptan sodium phosphate.
Notes: Drugs with a new active ingredient indicated for the treatment of fluid retention in heart failure patients who have not responded sufficiently to other diuretics to such as loop diuretics.